PMDA approval: Actonel Tablets 75 mg (Ajinomoto Pharmaceutical Co., Ltd.) Benet Tablets 75 mg
Applicant: Takeda Pharmaceutical Company Limited. Approval date: 2012-12-25. Type: New approval. Active ingredient(s): Sodium risedronate hydrate.
Notes: Drugs with a new dosage in a new additional dosage form. These drugs are indicated for the treatment of osteoporosis.